PMDA approval: Eliquis Tablets 2.5 mg Eliquis Tablets 5 mg
Applicant: Bristol-Myers K.K. Approval date: 2015-12-21. Type: Partial change. Active ingredient(s): Apixaban.
Notes: Drugs with a new additional indication and a new dosage for the treatment and prevention of the recurrence of venous thromboembolism (deep vein thrombosis and pulmonary thromboembolism).